PMDA approval: OneDuro Patch 0.84 mg OneDuro Patch 1.7 mg OneDuro Patch 3.4 mg OneDuro Patch 5 mg OneDuro Patch 6.7 mg
Applicant: Janssen Pharmaceutical K.K. Approval date: 2010-10-27. Type: New approval. Active ingredient(s): Fentanyl.
Notes: Drugs in new dosage forms indicated for the analgesia in various types of cancer with moderate to severe pain which cannot be managed by treatments with non-opioid analgesics and weak opioid analgesics (for use only in patients who switch from an opioid analgesic).